PMDA approval: Cravit Intravenous Drip Infusion 500 mg/100 mL Cravit Intravenous Drip Infusion 500 mg/20 mL
Applicant: Daiichi Sankyo Company, Limited. Approval date: 2010-10-27. Type: New approval. Active ingredient(s): Levofloxacin hydrate.
Notes: Drugs with a new route of administration indicated for the treatment of pneumonia, secondary infection of chronic respiratory disease, typhoid, paratyphoid, anthrax, brucellosis, plague, tularemia and Q fever.